PMDA approval: Nurtec OD Tablets 75 mg
Applicant: Pfizer Japan Inc. Approval date: 2025-09-19. Type: New approval. Active ingredient(s): Rimegepant sulfate hydrate.
Notes: A drug with a new active ingredient indicated for the acute treatment and prevention of migraine attacks.